PMDA approval: Remicade for I.V. Infusion 100
Applicant: Mitsubishi Tanabe Pharma Corporation. Approval date: 2011-08-17. Type: Partial change. Active ingredient(s): Infliximab (genetical recombination).
Notes: A drug with a new dosage indicated for the treatment of Crohn's disease. [Orphan drug]